PMDA approval: Trintellix Tablets 10 mg Trintellix Tablets 20 mg
Applicant: Takeda Pharmaceutical Company Limited. Approval date: 2019-09-20. Type: New approval. Active ingredient(s): Vortioxetine hydrobromide.
Notes: Drugs with a new active ingredient indicated for the treatment of depression.